PMDA approval: Forxiga 5 mg Tablets Forxiga 10 mg Tablets
Applicant: AstraZeneca K.K. Approval date: 2019-03-26. Type: Partial change. Active ingredient(s): Dapagliflozin propylene glycolate hydrate.
Notes: Drugs with a new additional indication and a new dosage for the treatment of type 1 diabetes mellitus.